PMDA approval: Tasfygo Tablets 35 mg
Applicant: Eisai Co., Ltd. Approval date: 2024-09-24. Type: New approval. Active ingredient(s): Tasurgratinib succinate.
Notes: A drug with a new active ingredient indicated for the treatment of unresectable FGFR2 fusion gene-positive biliary tract cancer that has progressed after cancer chemotherapy. [Orphan drug]